PMDA approval: Endoxan Tablets 50mg
Applicant: Shionogi & Co., Ltd. Approval date: 2022-02-25. Type: Partial change. Active ingredient(s): Cyclophosphamide hydrate.
Notes: A drug with other characteristics indicated for the suppression of immunoreaction associated with cell transplantation. [Expedited review]